PMDA approval: Rixubis Intravenous 250 Rixubis Intravenous 500 Rixubis Intravenous 1000 Rixubis Intravenous 2000 Rixubis Intravenous 3000
Applicant: Baxter Limited. Approval date: 2014-12-26. Type: New approval. Active ingredient(s): Nonacog gamma (genetical recombination).
Notes: Drugs with a new active ingredient indicated for the control of bleeding tendency in patients with blood coagulation factor IX deficiency.